PMDA approval: Botox for injection 50 Units Botox for Injection 100 Units
Applicant: GlaxoSmithKline K.K. Approval date: 2015-06-26. Type: Partial change. Active ingredient(s): Botulinum toxin type A.
Notes: Drugs with a new additional indication and a new dosage for the treatment of strabismus.